PMDA approval: Idelvion I.V. Injection 250 Idelvion I.V. Injection 500 Idelvion I.V. Injection 1000 Idelvion I.V. Injection 2000
Applicant: CSL Behring K.K. Approval date: 2016-09-28. Type: New approval. Active ingredient(s): Albutrepenonacog alfa (genetical recombination).
Notes: Drugs with a new active ingredient indicated for the control of bleeding tendency in patients with coagulation factor IX deficiency.